PMDA approval: Growject BC for Injection 8 mg Growject for Injection 8 mg Growject for Injection 1.33 mg
Applicant: JCR Pharmaceuticals Co., Ltd. Approval date: 2012-08-24. Type: Partial change. Active ingredient(s): Somatropin (genetical recombination).
Notes: Drugs with a new additional indication and a new dosage for the treatment of SGA (small-for-gestational age) dwarfism not associated with epiphyseal closure.